PMDA approval: Voydeya Tablets 50 mg
Applicant: Alexion Pharma GK. Approval date: 2024-01-18. Type: New approval. Active ingredient(s): Danicopan.
Notes: A drug with a new active ingredient indicated for the treatment of paroxysmal nocturnal haemoglobinuria. [Orphan drug]